PMDA approval: Cyramza Injection 100 mg Cyramza Injection 500 mg
Applicant: Eli Lilly Japan K.K. Approval date: 2015-03-26. Type: New approval. Active ingredient(s): Ramucirumab (genetical recombination).
Notes: Drugs with a new active ingredient indicated for the treatment of unresectable advanced or recurrent gastric cancer. [Priority review]